PMDA approval: Lasvic Intravenous Drip Infusion Kit 150 mg
Applicant: Kyorin Pharmaceutical Co., Ltd. Approval date: 2020-11-27. Type: New approval. Active ingredient(s): Lascufloxacin hydrochloride.
Notes: A drug with a new route of administration indicated for the treatment of pneumonia, lung abscess, and secondary infection of chronic respiratory disease caused by Lasvic-sensitive Staphylococcus spp, Streptococcus spp, Streptococcus pneumoniae, Enterococcus spp, Moraxella (Branhamella ) catarrhalis, Escherichia coli, Klebsiella spp, Enterobacter spp, Haemophilus influenzae, Legionella pneumophila, Peptostreptococcus spp, Veillonella spp, Bacteroides spp, Prevotella spp, Porphyromonas spp, Fusobacterium spp, and Mycoplasma pneumoniae.